PMDA approval: Botox for Injection 50 Units Botox for Injection 100 Units
Applicant: GlaxoSmithKline K.K. Approval date: 2024-06-24. Type: Partial change. Active ingredient(s): Botulinum toxin type A.
Notes: Drugs with a new indication and a new additional pediatric dosage for the treatment of upper and lower limb spasticity.